PMDA approval: Zykadia Capsules 150 mg
Applicant: Novartis Pharma K.K. Approval date: 2019-02-21. Type: Partial change. Active ingredient(s): Ceritinib.
Notes: A drug with a new dosage indicated for the treatment of unresectable advanced/relapsed ALK fusion gene-positive non-small-cell lung cancer.